PMDA approval: Anti-D Human Immunoglobulin I.M. 1000-Benesis
Applicant: Benesis Corporation. Approval date: 2011-05-20. Type: Partial change. Active ingredient(s): Dry anti-Rho(D) immune human globulin.
Notes: A drug with a new indication and a new dosage for the prevention of sensitization to the D(Rho) factor in women who are D(Rho) negative and are not already sensitized to the D(Rho) factor, to be used at around 28 weeks of pregnancy or in the case of possible sensitization such as after delivery, miscarriage, artificial abortion, ectopic pregnancy, testing/procedure during pregnancy (e.g., amniocentesis, external cephalic version) or abdominal bruise. [Public knowledge-based application after PAFSC's preliminary assessment]